PMDA approval: Botox for Injection 50 Units Botox for Injection 100 Units
Applicant: GlaxoSmithKline K.K. Approval date: 2018-05-25. Type: Partial change. Active ingredient(s): Botulinum toxin type A.
Notes: Drugs with a new additional indication and a new dosage for the treatment of spasmodic dysphonia.